PMDA approval: Elplat I.V. Infusion Solution 50 mg Elplat I.V. Infusion Solution 100 mg Elplat I.V. Infusion Solution 200 mg
Applicant: Yakult Honsha Co., Ltd. Approval date: 2013-12-20. Type: Partial change. Active ingredient(s): Oxaliplatin.
Notes: Drugs with a new additional indication and a new dosage for the treatment of unresectable pancreatic cancer. [Priority review], [Expedited review]